PMDA approval: Filgrastim BS Inj.75 mg Syringe “Sandoz” Filgrastim BS Inj.150 mg Syringe “Sandoz” Filgrastim BS Inj.300 mg Syringe “Sandoz”
Applicant: Sandoz K.K. Approval date: 2014-03-24. Type: New approval. Active ingredient(s): Filgrastim (genetical recombination) [Filgrastim biosimilar 3].
Notes: Follow-on biologics indicated for mobilization of hematopoietic stem cell to peripheral blood, promotion of increase in neutrophil count at the time of hematopoietic stem cell transplantation, and the treatment of neutropenia caused by cancer chemotherapy, neutropenia which affects the treatment of human immunodeficiency virus (HIV) infection, neutropenia associated with myelodysplastic syndrome, neutropenia associated with aplastic anemia and congenital/idiopathic neutropenia.